PMDA approval: Ultiva Intravenous 2 mg Ultiva Intravenous 5 mg
Applicant: Janssen Pharmaceutical K.K. Approval date: 2016-08-26. Type: Partial change. Active ingredient(s): Remifentanil hydrochloride.
Notes: Drugs with a new additional indication and a new dosage for use in pediatric patients for analgesia during maintenance of general anesthesia.